PMDA approval: L-Cartin FF Oral Solution 10% L-Cartin FF Injection 1000 mg
Applicant: Otsuka Pharmaceutical Co., Ltd. Approval date: 2012-12-25. Type: New approval. Active ingredient(s): Levocarnitine.
Notes: Drugs with a new active ingredient indicated for the treatment of carnitine deficiency.